PMDA approval: FerriSeltz Powder 20%
Applicant: Otsuka Pharmaceutical Co., Ltd. Approval date: 2010-03-12. Type: Partial change. Active ingredient(s): Ferric ammonium citrate.
Notes: A drug with a new additional indication and a new dosage for negative contrast of digestive tract in cholangiopancreatography.